PMDA approval: Oncovin for Inj. 1 mg
Applicant: Nippon Kayaku Co., Ltd. Approval date: 2013-03-25. Type: Partial change. Active ingredient(s): Vincristine sulfate.
Notes: A drug with a new additional indication and a new dosage for the treatment of pheochromocytoma. [Public knowledge-based application after PAFSC's preliminary assessment]